PMDA approval: Tezspire Subcutaneous Injection 210 mg Syringe
Applicant: AstraZeneca K.K. Approval date: 2022-09-26. Type: New approval. Active ingredient(s): Tezepelumab (genetical recombination).
Notes: A drug with a new active ingredient indicated for the treatment of bronchial asthma (for use only in patients with severe or intractable bronchial asthma whose asthmatic responses are uncontrollable with conventional therapies).